PMDA approval: Rituxan Intravenous Infusion 100 mg Rituxan Intravenous Infusion 500 mg
Applicant: Zenyaku Kogyo Co., Ltd. Approval date: 2024-09-24. Type: Partial change. Active ingredient(s): Rituximab (genetical recombination).
Notes: Drugs with a new indication and a new dosage for the treatment of refractory nephrotic syndrome (for use steroid-resistant nephrotic syndrome). [Orphan drug]